PMDA approval: Choreazine Tablets 12.5 mg
Applicant: Alfresa Pharma Corporation. Approval date: 2012-12-25. Type: New approval. Active ingredient(s): Tetrabenazine.
Notes: A drug with a new active ingredient indicated for the treatment of chorea associated with Huntington's disease. [Orphan drug]